PMDA approval: Balversa Tablets 3 mg Balversa Tablets 4 mg Balversa Tablets 5 mg
Applicant: Janssen Pharmaceutical K.K. Approval date: 2024-12-27. Type: New approval. Active ingredient(s): Erdafitinib.
Notes: Drugs with a new active ingredient indicated for the treatment of unresectable urothelial carcinoma with FGFR 3 gene mutation or fusion gene that has progressed after cancer chemotherapy.